PMDA approval: Pasil Intravenous Drip Infusion 300 mg Pasil Intravenous Drip Infusion 500 mg Pasil Intravenous Drip Infusion 1000 mg (Toyama Chemical Co., Ltd.) Pazucross Injection 300 Pazucross Injection 500 Pazucross Injection 1000 mg
Applicant: Mitsubishi Tanabe Pharma Corporation. Approval date: 2010-07-23. Type: Partial change. Active ingredient(s): Pazufloxacin mesilate.
Notes: Drugs with new additional indications for the treatment of Streptococcus pneumoniae and sepsis and a new dosage in an additional dosage form.